PMDA approval: Suiny Tab. 100 mg
Applicant: Sanwa Kagaku Kenkyusho Co., Ltd. Approval date: 2015-12-21. Type: Partial change. Active ingredient(s): Anagliptin.
Notes: A drug with a revised indication for the treatment of type 2 diabetes mellitus.